PMDA approval: Olumiant Tablets 4 mg Olumiant Tablets 2 mg
Applicant: Eli Lilly Japan K.K. Approval date: 2020-12-25. Type: Partial change. Active ingredient(s): Baricitinib.
Notes: Drugs with a new indication for the treatment of atopic dermatitis in patients who have not responded sufficiently to conventional treatments.